PMDA approval: MyoMIBG-I123 Injection
Applicant: Fujifilm RI Pharma Co., Ltd. Approval date: 2011-05-20. Type: Partial change. Active ingredient(s): 3-iodobenzylguanidine (123I) injection.
Notes: A drug with a new additional indication and a new dosage for diagnosis of melanocytoma in tumor scintigraphy. [Public knowledge-based application after PAFSC's preliminary assessment]